PMDA approval: Suprecur Nasal Solution 0.15%
Applicant: Sanofi K.K. Approval date: 2022-02-25. Type: Partial change. Active ingredient(s): Buserelin acetate.
Notes: A drug with a new indication and a new dosage for the induction of final follicular maturation in assisted reproductive technology. [Public knowledge-based application]